PMDA approval: Reclast for i.v. infusion 5 mg
Applicant: Asahi Kasei Pharma Corporation. Approval date: 2016-09-28. Type: New approval. Active ingredient(s): Zoledronic acid hydrate.
Notes: A drug with a new indication and a new dosage in an additional dosage form indicated for the treatment of osteoporosis.